PMDA approval: Nuwiq for I.V. Injection 250 Nuwiq for I.V. Injection 500 Nuwiq for I.V. Injection 1000 Nuwiq for I.V. Injection 2000 Nuwiq for I.V. Injection 2500 Nuwiq for I.V. Injection 3000 Nuwiq for I.V. Injection 4000
Applicant: Fujimoto Pharmaceutical Corporation. Approval date: 2021-01-22. Type: New approval. Active ingredient(s): Simoctocog alfa (genetical recombination).
Notes: Drugs with a new active ingredient indicated for the control of bleeding tendency in patients with blood coagulation factor VIII deficiency.